PMDA approval: Bipresso Extended Release Tablets 50 mg Bipresso Extended Release Tablets 150 mg
Applicant: Astellas Pharma Inc. Approval date: 2017-07-03. Type: New approval. Active ingredient(s): Quetiapine fumarate.
Notes: Drugs with a new indication in new dosage form indicated for the improvement of depressive symptoms in patients with bipolar disorder.